PMDA approval: Topina Topina Tablets 50 mg TopinaTablets 100 mg
Applicant: Kyowa Hakko Kogyo Co., Ltd. Approval date: 2007-07-31. Type: New approval. Active ingredient(s): Topiramate.
Notes: Drugs containing a new active ingredient indicated for use as an adjunctive therapy with other antiepileptic drugs to treat partial seizures (including secondary generalized seizures) in patients with epilepsy not fully responding to other antiepileptic therapies.